PMDA approval: Botox for Injection 50 Units Botox for Injection 100 Units
Applicant: GlaxoSmithKline K.K. Approval date: 2012-11-21. Type: Partial change. Active ingredient(s): Botulinum toxin type A.
Notes: Drugs with a new route of administration and an additional indication for the treatment of severe primary axillary hyperhidrosis.